PMDA approval: Anaemetro Intravenous Infusion 500 mg
Applicant: Pfizer Japan Inc. Approval date: 2014-07-04. Type: New approval. Active ingredient(s): Metronidazole.
Notes: A drug with a new route of administration indicated for the treatment of different kinds of anaerobic bacterial infections, infectious enteritis, and amebic dysentery.